PMDA approval: Cabometyx Tablets 20 mg Cabometyx Tablets 60 mg
Applicant: Takeda Pharmaceutical Company Limited. Approval date: 2020-11-27. Type: Partial change. Active ingredient(s): Cabozantinib malate.
Notes: Drugs with a new indication for the treatment of unresectable hepatocellular carcinoma that has progressed after cancer chemotherapy.